PMDA approval: Soliris for Intravenous Infusion 300 mg
Applicant: Alexion Pharma G.K. Approval date: 2017-12-25. Type: Partial change. Active ingredient(s): Eculizumab (genetical recombination).
Notes: A drug with a new additional indication and a new dosage indicated for the treatment of generalized myasthenia gravis (for use only in patients whose symptoms cannot be sufficiently controlled with high-dose intravenous immunoglobulin or plasmapheresis). [Orphan drug]